PMDA approval: Belsomra Tablets 15 mg Belsomra Tablets 20 mg
Applicant: MSD K.K. Approval date: 2014-09-26. Type: New approval. Active ingredient(s): Suvorexant.
Notes: Drugs with a new active ingredient indicated for the treatment of insomnia.